PMDA approval: G-Lasta Subcutaneous Injection 3.6 mg
Applicant: Kyowa Kirin Co., Ltd. Approval date: 2024-05-17. Type: Partial change. Active ingredient(s): Pegfilgrastim (genetical recombination).
Notes: A drug with a new indication for the mobilization of hematopoietic stem cells to the peripheral blood for autologous hematopoietic stem cell transplantation.